PMDA approval: Remicade for I.V. Infusion 100
Applicant: Mitsubishi Tanabe Pharma Corporation. Approval date: 2016-05-13. Type: Partial change. Active ingredient(s): Infliximab (genetical recombination).
Notes: A drug with a new dosage indicated for plaque psoriasis, psoriatic arthritis, pustular psoriasis, and erythrodermic psoriasis in patients who have not responded sufficiently to conventional treatments.